PMDA approval: Portrazza Injection 800 mg
Applicant: Eli Lilly Japan K.K. Approval date: 2019-06-18. Type: New approval. Active ingredient(s): Necitumumab (genetical recombination).
Notes: A drug with a new active ingredient indicated for the treatment of unresectable advanced or recurrent squamous non-small cell lung cancer.